PMDA approval: Spinraza Intrathecal Injection 28 mg Spinraza Intrathecal Injection 50 mg
Applicant: Biogen Japan Ltd. Approval date: 2025-09-19. Type: New approval. Active ingredient(s): Nusinersen sodium.
Notes: Drugs with a new dosage in an additional dosage form indicated for the treatment of spinal muscular atrophy. [Orphan drug]